PMDA approval: Endoxan for Injection 100 mg Endoxan for Injection 500 mg
Applicant: Shionogi & Co., Ltd. Approval date: 2013-03-25. Type: Partial change. Active ingredient(s): Cyclophosphamide hydrate.
Notes: Drugs with a new additional indication and a new dosage for the treatment of pheochromocytoma. [Public knowledge-based application after PAFSC's preliminary assessment]